PMDA approval: Lora-Pita Intravenous Injection 2 mg
Applicant: Pfizer Japan Inc. Approval date: 2018-09-21. Type: New approval. Active ingredient(s): Lorazepam.
Notes: A drug with a new route of administration indicated for the treatment of status epilepticus.